PMDA approval: Pariet Tablets 10 mg
Applicant: Eisai Co., Ltd. Approval date: 2010-06-18. Type: Partial change. Active ingredient(s): Sodium rabeprazole.
Notes: A drug with a new additional indication and a new dosage for the treatment of non-erosive reflux disease.